PMDA approval: Ondansetron Injection 4 mg Syringe “Maruishi”
Applicant: Maruishi Pharmaceutical Co., Ltd. Approval date: 2022-02-25. Type: Partial change. Active ingredient(s): Ondansetron hydrochloride hydrate.
Notes: A drug with a new indication and a new dosage for the treatment of digestive symptoms (nausea and vomiting) after surgery. [Public knowledge-based application after PAFSC's preliminary assessment]